PMDA approval: Kenketu Glovenin-I for I.V. Injection 500 mg Kenketu Glovenin-I for I.V. Injection 2500 mg Kenketu Glovenin-I for I.V. Injection 5000 mg
Applicant: Nihon Pharmaceutical Co., Ltd. Approval date: 2016-09-28. Type: Partial change. Active ingredient(s): Freeze-dried polyethylene glycol treated human normal immunoglobulin.
Notes: Drugs with a new additional indication for the treatment of Guillain-Barré syndrome (severe cases in an acute exacerbation phase with difficulty in walking).